PMDA approval: Lunabell Tablets
Applicant: Nobelpharma Co., Ltd. Approval date: 2008-04-16. Type: New approval. Active ingredient(s): Norethisterone/ ethinylestradiol.
Notes: A new combination drug indicated for the treatment of dysmenorrhea associated with endometriosis.